PMDA approval: Artcereb Irrigation and Perfusion Solution for Cerebrospinal Surgery
Applicant: Ostuka Pharmaceutical Factory, Inc. Approval date: 2007-10-19. Type: New approval. Active ingredient(s): N/A because of a new combination drug.
Notes: A new combination drug indicated for irrigation during craniotomy/trepanation and spinal surgery, and perfusion during endoscopic neurosurgery.